PMDA approval: Perjeta Intravenous Infusion 420 mg/14 mL
Applicant: Chugai Pharmaceutical Co., Ltd. Approval date: 2013-06-28. Type: New approval. Active ingredient(s): Pertuzumab (genetical recombination).
Notes: A drug with a new active ingredient indicated for the treatment of unresectable or recurrent HER2-positive breast cancer.